PMDA approval: Evrenzo Tablets 20 mg Evrenzo Tablets 50 mg Evrenzo Tablets 100 mg
Applicant: Astellas Pharma Inc. Approval date: 2020-11-27. Type: Partial change. Active ingredient(s): Roxadustat.
Notes: Drugs with a new indication for the treatment of renal anemia.